PMDA approval: Xeplion Aqueous Suspension for Intramuscular Injection 25 mg Syringe Xeplion Aqueous Suspension for Intramuscular Injection 50 mg Syringe Xeplion Aqueous Suspension for Intramuscular Injection 75 mg Syringe Xeplion Aqueous Suspension for Intramuscular Injection 100 mg Syringe Xeplion Aqueous Suspension for Intramuscular Injection 150 mg Syringe
Applicant: Janssen Pharmaceutical K.K. Approval date: 2013-09-20. Type: New approval. Active ingredient(s): Paliperidone palmitate.
Notes: Drugs with a new active ingredient indicated for the treatment of schizophrenia.